PMDA approval: Inderal Tablets 10 mg Inderal Tablets 20 mg
Applicant: AstraZeneca K.K. Approval date: 2014-11-18. Type: Partial change. Active ingredient(s): Propranolol hydrochloride.
Notes: Drugs with a new additional indication and a new dosage for the prevention of hypoxic attack caused by right ventricular outflow tract obstruction. [Public knowledge-based application after preliminary assessment by the Pharmaceutical Affairs and Food Sanitation Council (PAFSC)]